PMDA approval: Telavic 250 mg Tablet
Applicant: Mitsubishi Tanabe Pharma Corporation. Approval date: 2011-09-26. Type: New approval. Active ingredient(s): Telaprevir.
Notes: A drug with a new active ingredient indicated for the improvement of viremia in serogroup 1 (genotype I [1a] or II [1b]) chronic hepatitis C in: (1) untreated patients with high blood HCV RNA; or (2) patients who did not respond to, or in whom the symptom relapsed with, interferon monotherapy or interferon-ribavirin combination therapy. [Priority review]